PMDA approval: Emgality Subcutaneous Injection 120 mg Autoinjectors Emgality Subcutaneous Injection 120 mg Syringe
Applicant: Eli Lilly Japan K.K. Approval date: 2021-01-22. Type: New approval. Active ingredient(s): Galcanezumab (genetical recombination).
Notes: Drugs with a new active ingredient indicated for the prevention of migraine attacks.